PMDA approval: Tarceva Tablets 25 mg Tarceba Tablets 100 mg
Applicant: Chugai Pharmaceutical Co., Ltd. Approval date: 2011-07-01. Type: Partial change. Active ingredient(s): Erlotinib hydrochloride.
Notes: Drugs with a new additional indication and a new dosage for the treatment of unresectable pancreatic cancer.